PMDA approval: Rebetol Capsules 200 mg
Applicant: MSD K.K. Approval date: 2016-09-28. Type: Partial change. Active ingredient(s): Ribavirin.
Notes: A drug with a new additional indication and a new dosage for the improvement of viremia in patients with chronic hepatitis C in serogroup 2 (genotype 2), for a combination use of ombitasvir hydrate, paritaprevir hydrate, and ritonavir.